PMDA approval: Xeloda Tablets 300
Applicant: Chugai Pharmaceutical Co., Ltd. Approval date: 2007-12-12. Type: Partial change. Active ingredient(s): Capecitabine.
Notes: A drug with a new indication and dosage for use as a postoperative adjuvant chemotherapy of colon cancer. A drug with a new dosage for the treatment of inoperable or relapsed breast cancer.